PMDA approval: Estrana Tape 0.72 mg
Applicant: Hisamitsu Pharmaceutical Co., Inc. Approval date: 2014-02-21. Type: Partial change. Active ingredient(s): Estradiol.
Notes: A drug with a new additional indication and new dosages for the treatment of hypoestrogenism caused by hypogonadism, gonadectomy or primary ovarian insufficiency. [Public knowledge-based application after PAFSC's preliminary assessment]